PMDA approval: Velexbru Tablets 80 mg
Applicant: Ono Pharmaceutical Co., Ltd. Approval date: 2020-03-25. Type: New approval. Active ingredient(s): Tirabrutinib hydrochloride.
Notes: A drug with a new active ingredient indicated for the treatment of relapsed or refractory primary central nervous system (CNS) lymphoma. [Orphan drug]